PMDA approval: Buphenyl Tablet 500 mg Buphenyl Granule 94%
Applicant: CMIC Holdings, Co., Ltd. Approval date: 2012-09-28. Type: New approval. Active ingredient(s): Sodium phenylbutyrate.
Notes: Drugs with a new active ingredient indicated for the treatment of urea cycle disorder. [Orphan drug]